PMDA approval: Triumeq Combination Tablets
Applicant: ViiV Healthcare K.K. Approval date: 2015-03-16. Type: New approval. Active ingredient(s): Dolutegravir sodium, Abacavir sulfate, Lamivudine.
Notes: A new combination drug indicated for the treatment of HIV infection. [Orphan drug]